PMDA approval: Zerbaxa Combination for Intravenous Drip Infusion
Applicant: MSD K.K. Approval date: 2019-01-08. Type: New approval. Active ingredient(s): Ceftolozane sulfate/Tazobactam sodium.
Notes: A new combination drug with new active ingredients indicated for the treatment of cystitis, pyelonephritis, peritonitis, intra-abdominal abscess, cholecystitis, and liver abscess caused by Zerbaxa-sensitive Streptococcus spp., Escherichia coli, Citrobacter spp., Klebsiella spp., Enterobacter spp., Proteus spp., or Pseudomonas aeruginosa.